PMDA approval: Bi•Sifrol Tablets 0.125 mg Bi•Sifrol Tablets 0.5 mg
Applicant: Nippon Boehringer Ingelheim Co., Ltd. Approval date: 2010-01-20. Type: Partial change. Active ingredient(s): Pramipexole hydrochloride hydrate.
Notes: Drugs with a new additional indication and a new dosage for the treatment of moderate to severe idiopathic restless legs syndrome.